PMDA approval: Spevigo 450 mg for I.V. Infusion
Applicant: Nippon Boehringer Ingelheim Co., Ltd. Approval date: 2022-09-26. Type: New approval. Active ingredient(s): Spesolimab (genetical recombination).
Notes: A drug with a new active ingredient indicated for the improvement of acute symptoms in patients with pustular psoriasis.